PMDA approval: Lonsurf Combination Tablet T15 Lonsurf Combination Tablet T20
Applicant: Taiho Pharmaceutical Co., Ltd. Approval date: 2014-03-24. Type: New approval. Active ingredient(s): Trifluridine/tipiracil hydrochloride.
Notes: New combination drugs with a new active ingredient indicated for the treatment of unresectable advanced or recurrent colorectal cancer (for use only if refractory or intolerant to standard therapies).